PMDA approval: Thaled Capsule 100
Applicant: Fujimoto Pharmaceutical Corporation. Approval date: 2008-10-16. Type: New approval. Active ingredient(s): Thalidomide.
Notes: A drug containing a new active ingredient indicated for the treatment of relapsed or refractory multiple myeloma. [Orphan drug]